PMDA approval: Pentasa Tablets 250 mg Pentasa Tablets 500 mg
Applicant: Kyorin Pharmaceutical Co., Ltd. Approval date: 2012-08-24. Type: Partial change. Active ingredient(s): Mesalazine.
Notes: Drugs with a new dosage. These drugs are indicated for the treatment of ulcerative colitis.